PMDA approval: Yervoy Injection 50 mg
Applicant: Bristol-Myers Squibb K.K. Approval date: 2018-05-25. Type: Partial change. Active ingredient(s): Ipilimumab (genetical recombination).
Notes: A drug with a new dosage indicated for the treatment of unresectable melanoma. [Orphan drug]